PMDA approval: Actemra 80 mg for Intravenous Infusion Actemra 200 mg for Intravenous Infusion Actemra 400 mg for Intravenous Infusion
Applicant: Chugai Pharmaceutical Co., Ltd. Approval date: 2019-05-22. Type: Partial change. Active ingredient(s): Tocilizumab (genetical recombination).
Notes: Drugs with a new additional indication and a new dosage for the treatment of adult Still's disease in patients who have not responded sufficiently to conventional treatments.